PMDA approval: Jardiance Tablets 10 mg
Applicant: Nippon Boehringer Ingelheim Co., Ltd. Approval date: 2021-11-25. Type: Partial change. Active ingredient(s): Empagliflozin.
Notes: A drug with a new indication and a new dosage for the treatment of chronic heart failure (for use only in patients receiving standard treatment of chronic heart failure).